PMDA approval: Tenozet Tablets 300 mg
Applicant: GlaxoSmithKline K.K. Approval date: 2014-03-24. Type: New approval. Active ingredient(s): Tenofovir disoproxil fumarate.
Notes: A drug with a new active ingredient indicated for the growth inhibition of hepatitis B virus in patients with chronic hepatitis B who show liver dysfunction accompanied by proliferation of the virus. [Priority review]